PMDA approval: Movicol Combination Oral Solution
Applicant: EA Pharma Co., Ltd. Approval date: 2018-09-21. Type: New approval. Active ingredient(s): Macrogol 4000/Sodium chloride/Sodium bicarbonate/Potassium chloride.
Notes: A new combination drug indicated for the treatment of chronic constipation (excluding constipation due to organic diseases).